PMDA approval: Spiriva 2.5 μg Respimat 60 puffs Spiriva1.25 μg Respimat 60 puffs
Applicant: Nippon Boehringer Ingelheim Co., Ltd. Approval date: 2016-08-26. Type: Partial change. Active ingredient(s): Tiotropium bromide hydrate.
Notes: Drugs with a new additional indication, dosage, and dosage form for the relief of symptoms secondary to airway obstructive disorders in mild to moderate persistent bronchial asthma.